PMDA approval: Bimzelx Syringe for S.C Injection 160 mg Bimzelx Autoinjector for S.C Injection 160 mg
Applicant: UCB Japan Co., Ltd. Approval date: 2022-01-20. Type: New approval. Active ingredient(s): Bimekizumab (genetical recombination).
Notes: Drugs with a new active ingredient indicated for the treatment of plaque psoriasis, pustular psoriasis, and erythrodermic psoriasis in patients who have not responded sufficiently to conventional therapies.